PMDA approval: Kevzara 150 mg Syringe for SC Injection Kevzara 200 mg Syringe for SC Injection Kevzara 150 mg Auto-injector for SC Injection Kevzara 200 mg Auto-injector for SC Injection
Applicant: Sanofi K.K. Approval date: 2017-09-27. Type: New approval. Active ingredient(s): Sarilumab (genetical recombination).
Notes: Drugs with a new active ingredient indicated for the treatment of rheumatoid arthritis in patients who have not responded sufficiently to conventional treatments.